PMDA approval: Comirnaty RTU Intramuscular Injection
Applicant: Pfizer Japan Inc. Approval date: 2022-09-12. Type: Partial change. Active ingredient(s): Tozinameran Tozinameran and Riltozinameran.
Notes: A drug with a new indication, a new dosage, and other characteristics for the prevention of disease caused by SARS-CoV-2 infection (COVID-19). [Drug pertaining to Special Approval for Emergency]